PMDA approval: Abraxane I.V. Infusion 100 mg
Applicant: Taiho Pharmaceutical Co., Ltd. Approval date: 2010-07-23. Type: New approval. Active ingredient(s): Paclitaxel.
Notes: A drug in a new dosage form and with a new dosage indicated for the treatment of breast cancer.